PMDA approval: Spinraza Intrathecal Injection 12 mg
Applicant: Biogen Japan Ltd. Approval date: 2022-03-28. Type: Partial change. Active ingredient(s): Nusinersen sodium.
Notes: A drug with a new indication for the treatment of spinal muscular atrophy without any clinical manifestations that is predicted to develop with genetic test. [Orphan drug]